PMDA approval: Renail Gel 10%
Applicant: Maruho Co., Ltd. Approval date: 2023-03-27. Type: New approval. Active ingredient(s): Acetylcysteine.
Notes: A drug with a new route of administration indicated for the support of pincer nail correction.